PMDA approval: Soliris for Intravenous Infusion 300 mg
Applicant: Alexion Pharma GK. Approval date: 2023-08-23. Type: Partial change. Active ingredient(s): Eculizumab (genetical recombination).
Notes: A drug with a new additional pediatric dosage indicated for the treatment of generalized myasthenia gravis (for use only in patients whose symptoms cannot be sufficiently controlled with high-dose intravenous immunoglobulin therapy or plasmapheresis). [Orphan drug]